PMDA approval: Adlumiz Tablets 50 mg
Applicant: Ono Pharmaceutical Co., Ltd. Approval date: 2021-01-22. Type: New approval. Active ingredient(s): Anamorelin hydrochloride.
Notes: A drug with a new active ingredient indicated for the treatment of cancer cachexia in the following malignancies: non-small cell lung cancer, gastric cancer, pancreatic cancer, and colorectal cancer.